PMDA approval: Adsorbed Influenza Vaccine (H5N1) "Seiken" 1 mL
Applicant: Denka Seiken Co., Ltd. Approval date: 2013-03-25. Type: New approval. Active ingredient(s): Adsorbed influenza vaccine (H5N1).
Notes: A drug with a new active ingredient indicated for the prevention of pandemic influenza (H5N1). [Orphan drug]